PMDA approval: Nitopro Continuous Intravenous Solution 6 mg Nitopro Continuous Intravenous Solution 30 mg
Applicant: Maruishi Pharmaceutical Co., Ltd. Approval date: 2021-08-25. Type: Partial change. Active ingredient(s): Sodium nitroprusside hydrate.
Notes: Drugs with a new indication and a new dosage for the treatment of acute heart failure (including chronic heart failure in the exacerbation period) and hypertensive emergencies. [Public knowledge-based application after PAFSC's preliminary assessment]